PMDA approval: Zanosar for Intravenous Injection 1 g
Applicant: Nobelpharma Co., Ltd. Approval date: 2014-09-26. Type: New approval. Active ingredient(s): Streptozocin.
Notes: A drug with a new active ingredient indicated for the treatment of neuroendocrine tumors of the pancreas and gastrointestinal tract. [Orphan drug]